PMDA approval: Ranmark Subcutaneous Injection 120 mg
Applicant: Daiichi Sankyo Company, Limited. Approval date: 2012-01-18. Type: New approval. Active ingredient(s): Denosumab (genetical recombination).
Notes: A drug with a new active ingredient indicated for the treatment of bone lesions due to multiple myeloma and bone lesions due to bone metastasis of solid tumor.